PMDA approval: Alyssa Combination Tablets
Applicant: Fuji Pharma Co., Ltd. Approval date: 2024-09-24. Type: New approval. Active ingredient(s): Estetrol hydrate/Drospirenone.
Notes: A new combination drug with a new active ingredient indicated for the treatment of dysmenorrhea.